PMDA approval: Ninlaro Capsules 2.3 mg Ninlaro Capsules 3 mg Ninlaro Capsules 4 mg
Applicant: Takeda Pharmaceutical Company Limited. Approval date: 2020-03-25. Type: Partial change. Active ingredient(s): Ixazomib citrate.
Notes: Drugs with a new indication and a new dosage for the maintenance treatment of multiple myeloma in patients after autologous hematopoietic stem-cell transplantation.